PMDA approval: Gemzar Injection 200 mg Gemzar Injection 1 g
Applicant: Eli Lilly Japan K.K. Approval date: 2019-06-18. Type: Partial change. Active ingredient(s): Gemcitabine hydrochloride.
Notes: Drugs with a new dosage indicated for the treatment of non-small cell lung cancer.